PMDA approval: Hebsbulin-IH for Intravenous
Applicant: Benesis Corporation. Approval date: 2008-02-29. Type: Partial change. Active ingredient(s): Polyethylene glycol-treated human anti-HBs immunoglobulin.
Notes: A drug with a new indication and dosage for the prevention of hepatitis B recurrence in HBs antigen-positive recipients after liver transplantation and of hepatitis B occurrence in recipients after liver transplantation from HBc antigen-positive donors.